PMDA approval: (1) Olumiant Tablets 2 mg (2) Olumiant Tablets 4 mg (3) Olumiant Tablets 1 mg
Applicant: Eli Lilly Japan K.K. Approval date: 2024-03-26. Type: Partial change. Active ingredient(s): Baricitinib (1), (2).
Notes: Drugs with a new indication and a new dosage and (3) a drug with a new indication and a new dosage in an additional dosage form, for the treatment of polyarticular-course juvenile idiopathic arthritis in patients who have not responded sufficiently to conventional treatments.